PMDA approval: Carboplatin Injection 50 mg "NK" Carboplatin Injection 150 mg "NK" Carboplatin Injection 450 mg "NK"
Applicant: Mylan Seiyaku Ltd. Approval date: 2022-09-26. Type: Partial change. Active ingredient(s): Carboplatin.
Notes: Drugs with a new dosage indicated for the treatment of breast cancer.